PMDA approval: Regroth Dental Kit 600 μg Regroth Dental Kit 1200 μg
Applicant: Kaken Pharmaceutical Co., Ltd. Approval date: 2016-09-28. Type: New approval. Active ingredient(s): Trafermin (genetical recombination).
Notes: Drugs with a new route of administration indicated for the treatment of alveolar bone loss due to periodontitis.